PMDA approval: Eklira 400 μg Genuair 30 doses Eklira 400 μg Genuair 60 doses
Applicant: Kyorin Pharmaceutical Co., Ltd. Approval date: 2015-03-26. Type: New approval. Active ingredient(s): Aclidinium bromide.
Notes: Drugs with a new active ingredient indicated for the relief of symptoms secondary to airway obstructive disorder in chronic obstructive pulmonary disease (chronic bronchitis, emphysema).